PMDA approval: Rocephin Intravenous 0.5 g Rocephin Intravenous 1 g Rocephin Intravenous Drip 1 g Bag
Applicant: Chugai Pharmaceutical Co., Ltd. Approval date: 2007-11-13. Type: Partial change. Active ingredient(s): Ceftriaxone sodium.
Notes: Drugs with a new dosage for use in children, premature infants, and neonates.